PMDA approval: Cellcept Capsule 250
Applicant: Chugai Pharmaceutical Co., Ltd. Approval date: 2011-09-16. Type: Partial change. Active ingredient(s): Mycophenolate mofetil.
Notes: A drug with a new additional pediatric dosage indicated for the prevention of rejection in renal transplantation. [Public knowledge-based application after PAFSC's preliminary assessment]